PMDA approval: GalliaPharm 68Ge/68Ga Generator
Applicant: Eckert & Ziegler Radiopharma GmbH. Approval date: 2025-09-19. Type: New approval. Active ingredient(s): Gallium (68Ga) chloride.
Notes: A drug with a new active ingredient indicated for gallium (68Ga) radiolabelling of radiopharmaceutical precursors approved for positron emission tomography (PET) imaging.